PMDA approval: Lenvima Capsules 4 mg Lenvima Capsules 10 mg
Applicant: Eisai Co., Ltd. Approval date: 2021-12-24. Type: Partial change. Active ingredient(s): Lenvatinib mesilate.
Notes: Drugs with a new indication and a new dosage for the treatment of unresectable advanced or recurrent endometrial carcinoma that has progressed after cancer chemotherapy. [Orphan drug]